PMDA approval: Cosentyx for S.C. Injection 150 mg Syringe Cosentyx for S.C. Injection 150 mg Pen Cosentyx for S.C. Injection 75 mg Syringe
Applicant: Novartis Pharma K.K. Approval date: 2021-09-27. Type: Partial change. Active ingredient(s): Secukinumab (genetical recombination).
Notes: Drugs with a new additional pediatric dosage and a drug in an additional dosage form indicated for the treatment of plaque psoriasis, psoriatic arthritis, and pustular psoriasis in patients who have not responded sufficiently to conventional treatments.